PMDA approval: Taltz 80 mg Syringe for SC Injection Taltz 80 mg Auto-injector for SC Injection
Applicant: Eli Lilly Japan K.K. Approval date: 2020-09-25. Type: Partial change. Active ingredient(s): Ixekizumab (genetical recombination).
Notes: Drugs with a new indication and a new dosage for the treatment of non-radiographic axial spondyloarthritis in patients who have not responded sufficiently to conventional treatments.